PMDA approval: Ilaris for S.C. Injection 150 mg Ilaris Solution for S.C. Injection 150 mg
Applicant: Novartis Pharma K.K. Approval date: 2018-07-02. Type: Partial change. Active ingredient(s): Canakinumab (genetical recombination).
Notes: Drugs with a new additional indication and a new dosage for the treatment of systemic juvenile idiopathic arthritis in patients who have not responded sufficiently to conventional treatments. [Orphan drug]